PMDA approval: Zyprexa Tablets 2.5 mg Zyprexa Tablets 5 mg Zyprexa Tablets 10 mg Zyprexa Fine Granules 1% Zyprexa Zydis Tablets 5 mg Zyprexa Zydis Tablets 10 mg
Applicant: Eli Lilly Japan K.K. Approval date: 2010-10-27. Type: Partial change. Active ingredient(s): Olanzapine.
Notes: Drugs with a new additional indication and a new dosage for the improvement of manic symptoms in patients with bipolar disorder.